PMDA approval: Arzerra for I.V. infusion 100 mg Arzerra for I.V. infusion 1000 mg
Applicant: GlaxoSmithKline K.K. Approval date: 2013-03-25. Type: New approval. Active ingredient(s): Ofatumumab (genetical recombination).
Notes: Drugs with a new active ingredient indicated for the treatment of relapsed or refractory CD20-positive chronic lymphocytic leukemia. [Orphan drug]